PMDA approval: Calquence Tablets 100 mg
Applicant: AstraZeneca K.K. Approval date: 2025-08-25. Type: Partial change. Active ingredient(s): Acalabrutinib maleate hydrate.
Notes: A drug with a new indication and a new dosage for the treatment of mantle cell lymphoma.